PMDA approval: Hemangiol Syrup for Pediatric 0.375%
Applicant: Maruho Co., Ltd. Approval date: 2016-07-04. Type: New approval. Active ingredient(s): Propranolol hydrochloride.
Notes: A drug with a new indication in a new dosage form indicated for the treatment of infantile hemangioma. [Orphan drug]